PMDA approval: Maintate Tablets 2.5 Maintate Tablets 5 Maintate Tablets 0.625
Applicant: Mitsubishi Tanabe Pharma Corporation. Approval date: 2011-05-20. Type: Partial change. Active ingredient(s): Bisoprolol fumarate.
Notes: Drugs with a new additional indication and new dosages for the treatment of chronic heart failure secondary to ischemic heart disease or dilated cardiomyopathy in patients receiving basic treatment with angiotensin converting enzyme inhibitors or angiotensin II receptor antagonists, diuretics, digitalis preparations, etc. [Public knowledge-based application after PAFSC's preliminary assessment]